PMDA approval: Dinagest Tablets 0.5 mg
Applicant: Mochida Pharmaceutical Co., Ltd. Approval date: 2020-01-23. Type: New approval. Active ingredient(s): Dienogest.
Notes: A drug with a new indication and a new dosage in an additional dosage form for the treatment of dysmenorrhea.